PMDA approval: Foscarvir Infusion Solution 24 mg/mL
Applicant: AstraZeneca K.K. Approval date: 2011-05-20. Type: Partial change. Active ingredient(s): Foscarnet sodium hydrate.
Notes: A drug with new additional indications and a new dosage for the treatment of cytomegalovirus viremia and cytomegalovirus infection in patients undergoing hematopoietic stem cell transplantation.